PMDA approval: Humira 40 mg for S.C. Injection Syringe 0.8 mL
Applicant: Abbott Japan Co., Ltd. Approval date: 2010-01-20. Type: Partial change. Active ingredient(s): Adalimumab (genetical recombination).
Notes: A drug with new additional indications and a new dosage for the treatment of plaque psoriasis and psoriatic arthritis in patients who have not responded sufficiently to conventional treatments.